PMDA approval: Velcade Injection 3 mg
Applicant: Janssen Pharmaceutical K.K. Approval date: 2021-08-25. Type: Partial change. Active ingredient(s): Bortezomib.
Notes: A drug with a new indication and a new dosage for the treatment of systemic AL amyloidosis. [Orphan drug]